PMDA approval: Opfolda Capsules 65 mg
Applicant: Amicus Therapeutics, Inc. Approval date: 2025-06-24. Type: New approval. Active ingredient(s): Miglustat.
Notes: A drug with a new indication and a new dosage in an additional dosage form for use as a combination therapy with cipaglucosidase alfa (genetical recombination) for the treatment of late-onset Pompe disease (LOPD). [Orphan drug]